PMDA approval: Inovelon Tablets 100 mg Inovelon Tablets 200 mg
Applicant: Eisai Co., Ltd. Approval date: 2013-03-25. Type: New approval. Active ingredient(s): Rufinamide.
Notes: Drugs with a new active ingredient indicated for use as a concomitant therapy with other antiepileptic drugs to treat tonic and atonic seizures in patients with Lennox-Gastaut syndrome who have not responded sufficiently to other antiepileptic drugs. [Orphan drug]